PMDA approval: Mozobil Injection 24 mg
Applicant: Sanofi K.K. Approval date: 2016-12-19. Type: New approval. Active ingredient(s): Plerixafor.
Notes: A drug with a new active ingredient indicated for the enhancement of mobilization of hematopoietic stem cells to the peripheral blood for autologous peripheral blood stem cell transplantation. [Orphan drug]